PMDA approval: Liovel Combination Tablets LD Liovel Combination Tablets HD
Applicant: Takeda Pharmaceutical Company Limited. Approval date: 2011-07-01. Type: New approval. Active ingredient(s): Alogliptin benzoate/pioglitazone hydrochloride.
Notes: New combination drugs indicated for the treatment of type 2 diabetes mellitus.